PMDA approval: Keytruda Injection 100 mg
Applicant: MSD K.K. Approval date: 2022-08-24. Type: Partial change. Active ingredient(s): Pembrolizumab (genetical recombination).
Notes: A drug with a new indication and a new dosage for postoperative adjuvant treatment for renal cell carcinoma.